PMDA approval: Dinagest Tablets 1 mg Dinagest OD Tablets 1 mg
Applicant: Mochida Pharmaceutical Co., Ltd. Approval date: 2016-12-02. Type: Partial change. Active ingredient(s): Dienogest.
Notes: Drugs with a new additional indication for the alleviation of pain associated with uterine adenomyosis.